PMDA approval: Arokaris I.V. Infusion 235 mg
Applicant: Taiho Pharmaceutical Co., Ltd. Approval date: 2022-03-28. Type: New approval. Active ingredient(s): Fosnetupitant chloride hydrochloride.
Notes: A drug with a new active ingredient indicated for the treatment of digestive symptoms (nausea and vomiting, including delayed phase) resulting from the administration of antineoplastic agents (cisplatin, etc.).